PMDA approval: Trodelvy for Injection 200 mg
Applicant: Gilead Sciences, Inc. Approval date: 2024-09-24. Type: New approval. Active ingredient(s): Sacituzumab govitecan (genetical recombination).
Notes: A drug with a new active ingredient indicated for the treatment of hormone receptor-negative and human epidermal growth factor receptor 2 (HER2)-negative unresectable or recurrent breast cancer in patients who have previously been treated with chemotherapy. [Priority review]